PMDA approval: Fentanyl Injection 0.1 mg Sankyo Fentanyl Injection 0.25 mg Sankyo
Applicant: Daiichi Sankyo Propharma Co., Ltd. Approval date: 2007-08-23. Type: Partial change. Active ingredient(s): Fentanyl citrate.
Notes: Drugs with a new dosage for use in pediatric patients for inducing general anesthesia and for inducing analgesia during general anesthesia. Expedited review.